PMDA approval: NovoThirteen i.v.injection 2500
Applicant: Novo Nordisk Pharma Ltd. Approval date: 2015-03-26. Type: New approval. Active ingredient(s): Catridecacog (genetical recombination).
Notes: A drug with a new active ingredient indicated for the control of bleeding tendency in patients with congenital blood coagulation factor XIII A-subunit deficiency. [Orphan drug]